PMDA approval: Suprane Liquid for Inhalation
Applicant: Baxter Limited. Approval date: 2011-04-22. Type: New approval. Active ingredient(s): Desflurane.
Notes: A drug with a new active ingredient indicated for the maintenance of general anesthesia.